PMDA approval: Coralan Tablets 2.5 mg Coralan Tablets 5 mg Coralan Tablets 7.5 mg
Applicant: Ono Pharmaceutical Co., Ltd. Approval date: 2019-09-20. Type: New approval. Active ingredient(s): Ivabradine hydrochloride.
Notes: Drugs with a new active ingredient indicated for the treatment of chronic heart failure with sinus rhythm and a resting heart rate of 75 bpm or greater at baseline ( for use only in patients receiving standard treatment of chronic heart failure, including β-blocker).